PMDA approval: Risperdal Tablets 1 mg Risperdal Tablets 2 mg Risperdal Fine Granules 1% Risperdal OD Tablets 0.5 mg Risperdal OD Tablets 1 mg Risperdal OD Tablets 2 mg Risperdal Oral Solution 1 mg/mL
Applicant: Janssen Pharmaceutical K.K. Approval date: 2016-02-29. Type: Partial change. Active ingredient(s): Risperidone.
Notes: Drugs with a new additional indication and a new dosage for the treatment of irritability associated with autism spectrum disorder in children and adolescents.